PMDA approval: rHSA Bulk Solution Medway Injection 25% Medway Injection 5% (Mitsubishi Tanabe Pharma Corporation) rHSA Bulk Solution—Bipha Stem Injection 25% Stem Injection 5%
Applicant: Bipha Corporation. Approval date: 2007-10-19. Type: New approval. Active ingredient(s): Human serum albumin (genetical recombination).
Notes: Drugs containing a new active ingredient indicated for the treatment of hypoalbuminemia caused by loss of albumin (due to thermal burn, nephrotic syndrome, etc.) and reduced albumin synthesis (cirrhosis, etc.), and hemorrhagic shock.